PMDA approval: Amenalief Tablets 200 mg
Applicant: Maruho Co., Ltd. Approval date: 2023-02-24. Type: Partial change. Active ingredient(s): Amenamevir.
Notes: A drug with a new indication and a new dosage for the treatment of recurrent herpes simplex.